PMDA approval: Rituxan Injection 10 mg/mL
Applicant: Zenyaku Kogyo Co., Ltd. Approval date: 2016-02-29. Type: Partial change. Active ingredient(s): Rituximab (genetical recombination).
Notes: A drug with a new additional indication and a new dosage for prophylaxis of antibody-mediated rejection in patients who underwent ABO-incompatible kidney or liver transplantation. [Orphan drug]